PMDA approval: Valtrex Tablets 500 Valtrex Granules 50%
Applicant: GlaxoSmithKline K.K. Approval date: 2007-09-28. Type: Partial change. Active ingredient(s): Valaciclovir hydrochloride Valtrex Tablets 500.
Notes: A drug with a new indication and dosage for the treatment of varicella in adults and children.